PMDA approval: Velcade Injection 3 mg
Applicant: Janssen Pharmaceutical K.K. Approval date: 2019-08-22. Type: Partial change. Active ingredient(s): Bortezomib.
Notes: A drug with a new dosage indicated for the treatment of multiple myeloma. [Orphan drug]